PMDA approval: (1) Trulicity Subcutaneous Injection 0.75 mg Ateos (2) Trulicity Subcutaneous Injection 1.5 mg Ateos
Applicant: Eli Lilly Japan K.K. Approval date: 2024-06-24. Type: Partial change. Active ingredient(s): Dulaglutide (genetical recombination).
Notes: （1） A drug with a new dosage for the treatment of type 2 diabetes mellitus. （2） A drug with a new dosage in an additional dosage form for the treatment of type 2 diabetes mellitus.